PMDA approval: Zinplava for Intravenous Infusion 625 mg
Applicant: MSD K.K. Approval date: 2017-09-27. Type: New approval. Active ingredient(s): Bezlotoxumab (genetical recombination).
Notes: A drug with a new active ingredient indicated for the prevention of recurrent Clostridium difficile infection.